PMDA approval: Corebeta for Intravenous 12.5 mg
Applicant: Ono Pharmaceutical Co., Ltd. Approval date: 2011-07-01. Type: New approval. Active ingredient(s): Landiolol hydrochloride.
Notes: A drug with a new indication, a new dosage, and an additional dosage form indicated for the improvement of visualization of coronary arteries at high heart rate in coronary angiography by computed tomography.